PMDA approval: Opsumit Tablet 10 mg
Applicant: Actelion Pharmaceuticals Japan Ltd. Approval date: 2015-03-26. Type: New approval. Active ingredient(s): Macitentan.
Notes: A drug with a new active ingredient indicated for the treatment of pulmonary arterial hypertension.